PMDA approval: Januvia Tablets 12.5 mg Januvia Tablets 25 mg Januvia Tablets 50 mg Januvia Tablets 100 mg (MSD K.K.) Glactiv Tablets 12.5 mg Glactiv Tablets 25 mg Glactiv Tablets 50 mg Glactiv Tablets 100 mg
Applicant: Ono Pharmaceutical Co., Ltd. Approval date: 2014-05-23. Type: Partial change. Active ingredient(s): Sitagliptin phosphate hydrate.
Notes: Drugs with a new indication for the treatment of type 2 diabetes mellitus.